PMDA approval: Benlysta for S.C. Injection 200 mg Autoinjector
Applicant: GlaxoSmithKline K.K. Approval date: 2024-06-24. Type: Partial change. Active ingredient(s): Belimumab (genetical recombination).
Notes: A drug with a new additional pediatric dosage indicated for the treatment of systemic lupus erythematosus in patients who have not responded sufficiently to conventional treatments.